PMDA approval: Enhertu for Intravenous Drip Infusion 100 mg
Applicant: Daiichi Sankyo Company, Limited. Approval date: 2023-08-23. Type: Partial change. Active ingredient(s): Trastuzumab deruxtecan (genetical recombination).
Notes: A drug with a new indication for the treatment of unresectable advanced or recurrent HER2 (ERBB2 ) mutation-positive non-small cell lung cancer that has progressed after cancer chemotherapy. [Orphan drug]